PMDA approval: Atectura Inhalation Capsules Low Dose Atectura Inhalation Capsules Medium Dose Atectura Inhalation Capsules High Dose
Applicant: Novartis Pharma K.K. Approval date: 2020-06-29. Type: New approval. Active ingredient(s): Indacaterol acetate/Mometasone furoate.
Notes: New combination drugs with a new active ingredient indicated for the treatment of bronchial asthma (when a combination treatment of an inhaled steroid and a long-acting beta-2 agonist is needed).